PMDA approval: Ultomiris for Intravenous Infusion 300 mg Ultomiris for Intravenous Infusion 300 mg/3 mL Ultomiris for Intravenous Infusion 1100 mg/11 mL
Applicant: Alexion Pharma GK. Approval date: 2023-05-25. Type: Partial change. Active ingredient(s): Ravulizumab (genetical recombination).
Notes: Drugs with a new indication for the prevention of relapse of neuromyelitis optica spectrum disorder (including neuromyelitis optica).